PMDA approval: Aredia for I.V. Infusion 15 mg Aredia for I.V. Infusion 30 mg
Applicant: Novartis Pharma K.K. Approval date: 2014-05-23. Type: Partial change. Active ingredient(s): Pamidronate disodium hydrate.
Notes: Drugs with a new additional indication and a new dosage for the treatment of osteogenesis imperfecta. [Public knowledge-based application after PAFSC's preliminary assessment]